PMDA approval: Lixiana Tablets 15 mg Lixiana Tablets 30 mg Lixiana Tablets 60 mg Lixiana OD Tablets 15 mg Lixiana OD Tablets 30 mg Lixiana OD Tablets 60 mg
Applicant: Daiichi Sankyo Company, Limited. Approval date: 2025-02-20. Type: Partial change. Active ingredient(s): Edoxaban tosilate hydrate.
Notes: Drugs with a new indication for the prevention of thrombus/embolus formation in patients with chronic thromboembolic pulmonary hypertension.